PMDA approval: Riona Tab. 250 mg
Applicant: Japan Tobacco Inc. Approval date: 2014-01-17. Type: New approval. Active ingredient(s): Ferric citrate hydrate.
Notes: A drug with a new active ingredient indicated for the improvement of hyperphosphatemia in patients with chronic kidney disease.